PMDA approval: Rheumatrex Capsules 2 mg (Wyeth K.K.) Trexamette Capsules 2 mg (Shiono Chemical Co., Ltd.) Methotrexate Cap. 2 mg “Sawai” (Sawai Pharmaceutical Co., Ltd.) Methotrexate Capsules 2 mg “Towa” (Towa Pharmaceutical Co., Ltd.) Methotrexate Cap. 2 mg “Mylan” (Mylan Seiyaku Ltd.) Metolate Tablets 2 mg (Santen Pharmaceutical Co., Ltd.) Methotrexate Tablets 2 mg "Tanabe"
Applicant: Mitsubishi Tanabe Pharma Corporation. Approval date: 2008-09-24. Type: Partial change. Active ingredient(s): Methotrexate.
Notes: Drugs with a new indication and a new dosage for the treatment of juvenile idiopathic arthritis accompanied by articular symptoms. [Expedited review]